PMDA approval: Prevenar 20 Suspension Liquid for Injection
Applicant: Pfizer Japan Inc. Approval date: 2024-03-26. Type: New approval. Active ingredient(s): Pneumococcal polysaccharide conjugated to CRM197 carrier protein.
Notes: A drug with a new active ingredient indicated for the prophylaxis of invasive pneumococcal disease (serotypes 1, 3, 4, 5, 6A, 6B, 7F, 8, 9V, 10A, 11A, 12F, 14, 15B, 18C, 19A, 19F, 22F, 23F, and 33F) in children.